PMDA approval: Cervarix
Applicant: GlaxoSmithKline K.K. Approval date: 2009-10-16. Type: New approval. Active ingredient(s): HPV-16 L1 VLP and HPV-18 L1 VLP.
Notes: A drug with a new active ingredient indicated for prevention of cervical cancer (squamous-cell carcinoma and adenocarcinoma) and its precursor lesions (cervical intraepithelial neoplasia [CIN] 2 and 3) associated with human papillomavirus (HPV) types 16 and 18 infection. [Priority review]